PMDA approval: Intelence Tablets 100 mg
Applicant: Janssen Pharmaceutical K.K. Approval date: 2008-12-25. Type: New approval. Active ingredient(s): Etravirine.
Notes: A drug containing a new active ingredient indicated for the treatment of HIV-1 infection. [Orphan drug]